PMDA approval: Sprycel Tablets 20 mg Sprycel Tablets 50 mg
Applicant: Bristol-Myers K.K. Approval date: 2011-06-16. Type: Partial change. Active ingredient(s): Dasatinib hydrate.
Notes: Drugs with a new additional indication and a new dosage for the treatment of chronic myelocytic leukemia.